PMDA approval: Norlevo Tablets 0.75 mg
Applicant: Sosei Co., Ltd. Approval date: 2011-02-23. Type: New approval. Active ingredient(s): Levonorgestrel.
Notes: A drug with a new additional indication and a new dosage for emergency contraception.